PMDA approval: Corectim Ointment 0.25% Corectim Ointment 0.5%
Applicant: Japan Tobacco Inc. Approval date: 2021-03-23. Type: New approval. Active ingredient(s): Delgocitinib.
Notes: Drugs with a new additional pediatric dosage and a drug in an additional dosage form indicated for the treatment of atopic dermatitis.